PMDA approval: Silgard 9 Aqueous Suspension for Intramuscular Injection Syringes
Applicant: MSD K.K. Approval date: 2023-03-08. Type: Partial change. Active ingredient(s): Human papillomavirus L1 protein virus-like particles (types 6, 11, 16, 18, 31, 33, 45, 52, and 58).
Notes: A drug with a new dosage indicated for the prevention of the following diseases caused by infection with human papillomavirus types 6, 11, 16, 18, 31, 33, 45, 52, and 58: • Cervical cancer (squamous cell carcinoma and adenosquamous carcinoma) and its precursor lesions (cervical intraepithelial neoplasia [CIN] 1, 2, and 3, and adenocarcinoma in situ [AIS]) • Vulvar intraepithelial neoplasia (VIN) 1, 2, and 3, and vaginal intraepithelial noeplasia (VaIN) 1, 2, and 3 • Condyloma acuminatum [Expedited review]